PMDA approval: Flutiform 50 Aerosol 56 Puffs Flutiform 50 Aerosol 120 Puffs
Applicant: Kyorin Pharmaceutical Co., Ltd. Approval date: 2020-06-29. Type: Partial change. Active ingredient(s): Fluticasone propionate/Formoterol fumarate hydrate.
Notes: Drugs with a new additional pediatric dosage indicated for the treatment of bronchial asthma (when a combination treatment of an inhaled steroid and a long-acting beta-2 agonist is needed).